PMDA approval: Jyseleca Tablets 100 mg Jyseleca Tablets 200 mg
Applicant: Gilead Sciences K.K. Approval date: 2022-03-28. Type: Partial change. Active ingredient(s): Filgotinib maleate.
Notes: Drugs with a new indication and a new dosage for the treatment and the maintenance therapy of moderate to severe ulcerative colitis (for use only in patients who have not sufficiently responded to conventional treatments).